PMDA approval: Asmanex Twisthaler 100 μg 60 doses Asmanex Twisthaler 200 μg 60 doses
Applicant: Schering-Plough K.K. Approval date: 2009-07-07. Type: New approval. Active ingredient(s): Mometasone furoate.
Notes: Drugs with a new route of administration indicated for the treatment of bronchial asthma.